PMDA approval: (1) Feburic Tablet 10 mg (2) Feburic Tablet 20 mg (2) Feburic Tablet 40 mg
Applicant: Teijin Pharma Limited. Approval date: 2023-06-26. Type: Partial change. Active ingredient(s): Febuxostat.
Notes: (1) A drug with a new additional pediatric dosage and other characteristics indicated for the treatment of gout and hyperuricemia. (2) Drugs with a new additional pediatric dosage indicated for the treatment of gout and hyperuricemia.